PMDA approval: Cell Culture-derived Influenza Emulsion HA Vaccine H5N1 for Intramuscular Injection “Kaketsuken”
Applicant: Kaketsuken [The Chemo-Sero-Therapeutic Research Institute]. Approval date: 2014-03-24. Type: New approval. Active ingredient(s): Cell culture-derived influenza emulsion HA vaccine (H5N1).
Notes: A drug with a new active ingredient indicated for the prevention of pandemic influenza (H5N1). [Orphan drug]